PMDA approval: Symbicort Turbuhaler 30 doses Symbicort Turbuhaler 60 doses
Applicant: AstraZeneca K.K. Approval date: 2012-08-10. Type: Partial change. Active ingredient(s): Budesonide/formoterol fumarate hydrate.
Notes: Drugs with a new additional indication and a new dosage for the relief of symptoms of chronic obstructive pulmonary diseases (chronic bronchitis, emphysema).